PMDA approval: Divigel 1 mg
Applicant: Pola Pharma Inc. Approval date: 2007-07-31. Type: New approval. Active ingredient(s): Estradiol.
Notes: A drug in a new dosage form, or gel, indicated for the treatment of vasomotor symptoms (hot flushes and sweating) associated with menopausal disorders and ovarian deficiency symptoms.